PMDA approval: Gobik Aqueous Suspension Syringes
Applicant: The Research Foundation for Microbial Diseases of Osaka University. Approval date: 2023-03-27. Type: New approval. Active ingredient(s): Protective antigen of Bordetella pertussis Diphtheria toxoid Tetanus toxoid Inactivated poliovirus types 1-3 (Sabin strain) Haemophilus influenzae type b oligosaccharide - diphtheria CRM197 conjugate.
Notes: A new combination drug with a new active ingredient indicated for the prevention of pertussis, diphtheria, tetanus, acute poliomyelitis, and infections caused by Haemophilus influenzae type b.